PMDA approval: Entresto Granules for Pediatric 12.5 mg Entresto Granules for Pediatric 31.25 mg
Applicant: Novartis Pharma K.K. Approval date: 2024-03-26. Type: New approval. Active ingredient(s): Sacubitril valsartan sodium hydrate.
Notes: Drugs with a new indication and a new additional pediatric dosage in an additional dosage form for the treatment of chronic heart failure.